PMDA approval: Cymbalta Capsules 20 mg Cymbalta Capsules 30 mg
Applicant: Shionogi & Co., Ltd. Approval date: 2015-05-26. Type: Partial change. Active ingredient(s): Duloxetine hydrochloride.
Notes: Drugs with a new additional indication and a new dosage for the treatment of pain associated with fibromyalgia.